PMDA approval: Phesgo Combination for Subcutaneous Injection MA Phesgo Combination for Subcutaneous Injection IN
Applicant: Chugai Pharmaceutical Co., Ltd. Approval date: 2023-09-25. Type: New approval. Active ingredient(s): Pertuzumab (genetical recombination), Trastuzumab (genetical recombination), and Vorhyaluronidase alfa (genetical recombination).
Notes: New combination drugs with a new active ingredient indicated for the treatment of HER2-positive breast cancer and unresectable advanced or recurrent HER2-positive colon or rectal cancer that has progressed after cancer chemotherapy.